PMDA approval: Surepost Tablets 0.25 mg Surepost Tablets 0.5 mg
Applicant: Dainippon Sumitomo Pharma Co., Ltd. Approval date: 2013-02-28. Type: Partial change. Active ingredient(s): Repaglinide.
Notes: Drugs with a new indication for the improvement of postprandial changes of blood glucose in type 2 diabetes mellitus.